PMDA approval: Influenza HA Vaccine "Kaketsuken" TF Influenza HA Vaccine "Kaketsuken" Influ "Kaketsuken" Syringe
Applicant: Kaketsuken [The Chemo-Sero-Therapeutic Research Institute]. Approval date: 2011-08-08. Type: Partial change. Active ingredient(s): Influenza HA Vaccine.
Notes: Drugs with a new pediatric dosage indicated for the prevention of influenza. [Expedited review]